PMDA approval: MonoVer for I.V. Injection 500 mg MonoVer for I.V. Injection 1000 mg
Applicant: Nippon Shinyaku Co., Ltd. Approval date: 2022-03-28. Type: New approval. Active ingredient(s): Ferric derisomaltose.
Notes: Drugs with a new active ingredient indicated for the treatment of iron-deficiency anemia.